PMDA approval: Bicanate Infusion
Applicant: Ostuka Pharmaceutical Factory, Inc. Approval date: 2008-03-19. Type: New approval. Active ingredient(s): N/A because of a combination drug.
Notes: A combination drug similar to other products indicated for the supply/correction of extracellular fluid in the cases where circulating blood and interstitial fluid decrease, and for the correction of metabolic acidosis.